PMDA approval: Reminyl Tablets 4 mg Reminyl Tablets 8 mg Reminyl Tablets 12 mg Reminyl OD Tablets 4 mg Reminyl OD Tablets 8 mg Reminyl OD Tablets 12 mg Reminyl Oral Solution 4 mg/mL
Applicant: Janssen Pharmaceutical K.K. Approval date: 2011-01-21. Type: New approval. Active ingredient(s): Galantamine hydrobromide.
Notes: Drugs with a new active ingredient indicated for inhibition of progression of symptoms of dementia in mild and moderate Alzheimer's dementia.